PMDA approval: Tarceva Tablets 25 mg Tarceva Tablets 100 mg Tarceva Tablets 150 mg
Applicant: Chugai Pharmaceutical Co., Ltd. Approval date: 2007-10-19. Type: New approval. Active ingredient(s): Erlotinib.
Notes: Drugs containing a new active ingredient indicated for the treatment of unresectable, relapsed/advanced non-small cell lung cancer exacerbated after cancer chemotherapy. [Priority review]